PMDA approval: Cancidas for Intravenous Drip Infusion 50 mg Cancidas for Intravenous Drip Infusion 70 mg
Applicant: MSD K.K. Approval date: 2012-01-18. Type: New approval. Active ingredient(s): Caspofungin acetate.
Notes: Drugs with a new active ingredient indicated for the treatment of febrile neutropenia suspected of a fungal infection and fungal infections due to Candida or Aspergillus (esophageal candidiasis, invasive candidiasis, aspergillosis).